PMDA approval: Feiba NF for Injection 500 Feiba NF for Injection 1000
Applicant: Baxter Limited. Approval date: 2014-06-20. Type: Partial change. Active ingredient(s): Anti-inhibitor coagulant complex.
Notes: Drugs with a revised indication and a new dosage for inhibition of bleeding tendency by promoting the blood coagulation in the plasma in patients with inhibitors to blood coagulation factor VIII or factor IX.